PMDA approval: Diacomit Drysyrup 250 mg Diacomit Drysyrup 500 mg Diacomit Capsules 250 mg
Applicant: Meiji Seika Pharma Co., Ltd. Approval date: 2012-09-28. Type: New approval. Active ingredient(s): Stiripentol.
Notes: Drugs with a new active ingredient indicated for use in conjunction with clobazam and sodium valproate as adjunctive therapy of refractory generalized tonic-clonic and clonic seizures in patients with Dravet’s syndrome whose seizures are not adequately controlled with clobazam and sodium valproate. [Orphan drug]